PMDA approval: Vafseo Tablets 150 mg Vafseo Tablets 300 mg
Applicant: Mitsubishi Tanabe Pharma Corporation. Approval date: 2020-06-29. Type: New approval. Active ingredient(s): Vadadustat.
Notes: Drugs with a new active ingredient indicated for the treatment of renal anemia.